PMDA approval: Herceptin Intravenous Infusion 60 Herceptin Intravenous Infusion 150
Applicant: Chugai Pharmaceutical Co., Ltd. Approval date: 2022-03-28. Type: Partial change. Active ingredient(s): Trastuzumab (genetical recombination).
Notes: Drugs with a new indication and a new dosage for the treatment of unresectable advanced or recurrent HER2-positive colon or rectal cancer that has progressed after cancer chemotherapy.